PMDA approval: Prezcobix Combination Tablets
Applicant: Janssen Pharmaceutical K.K. Approval date: 2016-11-22. Type: New approval. Active ingredient(s): Darunavir ethanolate/ Cobicistat.
Notes: A new combination drug with a new active ingredient indicated for the treatment of HIV infection. [Orphan drug]